PMDA approval: Mikelan LA Ophthalmic Solution 1% Mikelan LA Ophthalmic Solution 2%
Applicant: Otsuka Pharmaceutical Co., Ltd. Approval date: 2007-04-18. Type: New approval. Active ingredient(s): Carteolol hydrochloride.
Notes: Drugs in a new dosage form indicated for the treatment of glaucoma and ocular hypertension.